PMDA approval: Rinvoq Tablets 15 mg Rinvoq Tablets 7.5 mg
Applicant: AbbVie GK. Approval date: 2025-06-24. Type: Partial change. Active ingredient(s): Upadacitinib hydrate.
Notes: Drugs with a new indication and a new dosage for the treatment of giant cell arteritis in patients who have not responded sufficiently to conventional treatments.